PMDA approval: Genvoya Combination Tablets
Applicant: Japan Tobacco Inc. Approval date: 2019-05-22. Type: Partial change. Active ingredient(s): Elvitegravir/Cobicistat /Emtricitabine/Tenofovir alafenamide fumarate.
Notes: A drug with a new additional pediatric dosage indicated for the treatment of HIV-1 infection. [Orphan drug]